PMDA approval: Concerta Tablets 18 mg Concerta Tablets 27 mg
Applicant: Janssen Pharmaceutical K.K. Approval date: 2007-10-26. Type: New approval. Active ingredient(s): Methylphenidate hydrochloride.
Notes: Drugs with a new indication for the treatment of attention-deficit/hyperactivity disorder (AD/HD) in children. Expedited review.